PMDA approval: 5-FU Injection 250 mg 5-FU Injection 1000 mg
Applicant: Kyowa Kirin Co., Ltd. Approval date: 2021-11-25. Type: Partial change. Active ingredient(s): Fluorouracil.
Notes: Drugs with a new indication and a new dosage for the treatment of esophageal cancer. [Public knowledge-based application]